PMDA approval: Nesina Tablets 6.25 mg Nesina Tablets 12.5 mg Nesina Tablets 25 mg
Applicant: Takeda Pharmaceutical Company Limited. Approval date: 2010-08-20. Type: Partial change. Active ingredient(s): Alogliptin benzoate.
Notes: Drugs with a new additional indication for the treatment of type 2 diabetes mellitus in patients who have not responded sufficiently to thiazolidinediones along with diet and exercise therapies.